PMDA approval: Typhim Vi Syringe for Injection
Applicant: Sanofi K.K. Approval date: 2024-06-24. Type: New approval. Active ingredient(s): Salmonella typhi (TY2 strain) purified Vi capsular polysaccharide.
Notes: A drug with a new active ingredient indicated for the prevention of typhoid fever.